PMDA approval: Adoair 100 Diskus Adoair 50 Air 120 puffs
Applicant: GlaxoSmithKline K.K. Approval date: 2009-01-21. Type: Partial change. Active ingredient(s): Salmeterol xinafoate/fluticasone propionate.
Notes: A drug with a new additional pediatric dosage in a new dosage form indicated for the treatment of bronchial asthma (for use when concomitant use of inhaled corticosteroid and long-acting β2-agonist is required). [Expedited review]